PMDA approval: Pegasys S.C. 180 μg Pegasys S.C. 90 μg
Applicant: Chugai Pharmaceutical Co., Ltd. Approval date: 2011-09-26. Type: Partial change. Active ingredient(s): Peginterferon alfa-2a (genetical recombination).
Notes: Drugs with a new additional indication and a new dosage for the improvement of viremia in patients with chronic active hepatitis B. [Priority review]